PMDA approval: Kaletra Combination Tablets
Applicant: Abbott Japan Co., Ltd. Approval date: 2010-12-08. Type: Partial change. Active ingredient(s): Lopinavir/ritonavir.
Notes: A drug with new dosages for the treatment of HIV infection. [Orphan drug]